PMDA approval: Paraplatin Injection 50 mg Paraplatin Injection 150 mg Paraplatin Injection 450 mg
Applicant: Clinigen K.K. Approval date: 2024-06-24. Type: Partial change. Active ingredient(s): Carboplatin.
Notes: Drugs with a new indication and a new dosage for the treatment of endometrial cancer. [Public knowledge-based application after PAFSC's preliminary assessment]